PMDA approval: Kerendia Tablets 10 mg Kerendia Tablets 20 mg
Applicant: Bayer Yakuhin, Ltd. Approval date: 2022-03-28. Type: New approval. Active ingredient(s): Finerenone.
Notes: Drugs with a new active ingredient indicated for the treatment of chronic kidney disease associated with type 2 diabetes mellitus (excluding patients who have end-stage renal failure or are undergoing dialysis).